PMDA approval: Amyvid Injection
Applicant: Fujifilm RI Pharma Co., Ltd. Approval date: 2016-12-19. Type: New approval. Active ingredient(s): Florbetapir (18F).
Notes: A drug with a new active ingredient indicated for visualization of beta ‑amyloid plaques in the brain of patients with cognitive impairment suspected to be Alzheimer's disease.